PMDA approval: (1) Briviact Tablets 25 mg (2) Briviact Tablets 50 mg (3) Briviact for I.V. Injection 25 mg
Applicant: UCB Japan Co., Ltd. Approval date: 2024-06-24. Type: New approval. Active ingredient(s): Brivaracetam (1) (2).
Notes: Drugs with a new active ingredient indicated for the treatment of partial seizures (including secondary generalized seizures) in patients with epilepsy. (3) A drug with a new active ingredient indicated for the following treatment; partial seizures (including secondary generalized seizures) in patients with epilepsy. It is used as an alternative therapy for blevaracetam oral formulation in patients who are temporarily unable to be administered orally.